PMDA approval: Humira 40 mg for S.C. Injection Syringe 0.8 mL Humira 40 mg for S.C. Injection Syringe 0.4 mL Humira 80 mg for S.C. Injection Syringe 0.8 mL
Applicant: AbbVie GK. Approval date: 2016-09-28. Type: Partial change. Active ingredient(s): Adalimumab (genetical recombination).
Notes: Drugs with a new additional indication and a new dosage for the treatment of non-infectious intermediate, posterior, and panuveitis in patients who have not responded sufficiently to conventional therapies.